PMDA approval: Rituxan Intravenous Infusion 100 mg Rituxan Intravenous Infusion 500 mg
Applicant: Zenyaku Kogyo Co., Ltd. Approval date: 2023-12-22. Type: Partial change. Active ingredient(s): Rituximab (genetical recombination).
Notes: Drugs with a new indication and a new dosage for the suppression of antibody-mediated rejection in the following organ transplantations : kidney transplantation, liver transplantation, heart transplantation, lung transplantation, pancreas transplantation, small intestine transplantation, and the treatment of antibody-mediated rejection in the following organ transplantations : kidney transplantation, liver transplantation, heart transplantation, lung transplantation, pancreas transplantation, small intestine transplantation. [Orphan drug]